PMDA approval: Bepio Gel 2.5%
Applicant: Maruho Co., Ltd. Approval date: 2014-12-26. Type: New approval. Active ingredient(s): Benzoyl peroxide.
Notes: A drug with a new active ingredient indicated for the treatment of acne vulgaris.